PMDA approval: Corectim Ointment 0.5%
Applicant: Japan Tobacco Inc. Approval date: 2020-01-23. Type: New approval. Active ingredient(s): Delgocitinib.
Notes: A drug with a new active ingredient indicated for the treatment of atopic dermatitis.